PMDA approval: Valtrex Tablets 500 Valtrex Granules 50%
Applicant: GlaxoSmithKline K.K. Approval date: 2014-11-18. Type: Partial change. Active ingredient(s): Valaciclovir hydrochloride.
Notes: Drugs with a new additional indication and revised indications with a new dosage for prevention of herpes simplex virus infection in adult or pediatric haematopoietic stem cell transplant patients, for treatment of herpes simplex /herpes zoster, and for prevention of recurrent genital herpes in pediatric patients.